PMDA approval: Remicade for I.V. Infusion 100
Applicant: Mitsubishi Tanabe Pharma Corporation. Approval date: 2015-08-24. Type: Partial change. Active ingredient(s): Infliximab (genetical recombination).
Notes: A drug with new additional indications and a new dosage for the treatment of entero-Behcet's disease, neuro-Behcet's disease, and vasculo-Behcet's disease in patients who have not responded sufficiently to conventional therapies. [Orphan drug]